PMDA approval: Somatuline Subcutaneous Injection 60 mg Somatuline Subcutaneous Injection 90 mg Somatuline Subcutaneous Injection 120 mg
Applicant: Teijin Pharma Limited. Approval date: 2012-06-29. Type: New approval. Active ingredient(s): Lanreotide acetate.
Notes: Drugs with a new active ingredient for the improvement of hypersecretion of growth hormone and IGF-I (somatomedin-C) and related symptoms in acromegaly and pituitary gigantism (when surgical therapies are not sufficiently effective or are difficult to perform).